PMDA approval: Leuplin for Injection 1.88 Leuplin for Injection 3.75
Applicant: Takeda Pharmaceutical Company Limited. Approval date: 2011-05-20. Type: Partial change. Active ingredient(s): Leuprorelin acetate.
Notes: Drugs with a new additional dosage indicated for the treatment of central precocious puberty. [Public knowledge-based application after PAFSC's preliminary assessment]